PMDA approval: Agalsidase Beta BS I.V. Infusion 5 mg [JCR] Agalsidase Beta BS I.V. Infusion 35 mg [JCR]
Applicant: JCR Pharmaceuticals Co., Ltd. Approval date: 2018-09-21. Type: New approval. Active ingredient(s): Agalsidase beta (genetical recombination) [agalsidase beta biosimilar 1].
Notes: Follow-on biologics indicated for the treatment of Fabry disease.